PMDA approval: Lonasen Tablets 2 mg Lonasen Tablets 4 mg Lonasen Powder 2%
Applicant: Dainippon Sumitomo Pharma Co., Ltd. Approval date: 2008-01-25. Type: New approval. Active ingredient(s): Blonanserin.
Notes: Drugs containing a new active ingredient indicated for the treatment of schizophrenia.